PMDA approval: 5-FU Injection 250 mg 5-FU Injection 1000 mg
Applicant: Kyowa Hakko Kirin Co., Ltd. Approval date: 2018-09-21. Type: Partial change. Active ingredient(s): Fluorouracil.
Notes: Drugs with a new indication and a new dosage for the treatment of small intestine cancer. [Public knowledge-based application after PAFSC's preliminary assessment]